PMDA approval: Verzenio Tablets 50 mg Verzenio Tablets 100 mg Verzenio Tablets 150 mg
Applicant: Eli Lilly Japan K.K. Approval date: 2018-09-21. Type: New approval. Active ingredient(s): Abemaciclib.
Notes: Drugs with a new active ingredient indicated for the treatment of hormone receptor (HR)-positive and human epidermal growth factor receptor 2 (HER2)-negative inoperable or recurrent breast cancer.